PMDA approval: Visiclear Tablets
Applicant: Zeria Pharmaceutical Co., Ltd. Approval date: 2007-04-18. Type: New approval. Active ingredient(s): Monobasic sodium phosphate monohydrate/dibasic sodium phosphate anhydrous.
Notes: A new combination drug indicated for bowel cleansing as a preparation for colonoscopy.